PMDA approval: Orladeyo Capsules 150 mg
Applicant: OrphanPacific, Inc. Approval date: 2021-01-22. Type: New approval. Active ingredient(s): Berotralstat hydrochloride.
Notes: A drug with a new active ingredient indicated for the prophylaxis of acute attacks of hereditary angioedema. [SAKIGAKE review, orphan drug]